PMDA approval: Nanozora 30 mg Syringes for S.C. Injection
Applicant: Taisho Pharmaceutical Co., Ltd. Approval date: 2022-09-26. Type: New approval. Active ingredient(s): Ozoralizumab (genetical recombination).
Notes: A drug with a new active ingredient indicated for the treatment of rheumatoid arthritis in patients who have not responded sufficiently to conventional treatments.